PMDA approval: Keytruda Injection 100 mg
Applicant: MSD K.K. Approval date: 2024-12-27. Type: Partial change. Active ingredient(s): Pembrolizumab (genetical recombination).
Notes: A drug with a new indication and a new dosage for the treatment of advanced or recurrent endometrial cancer. [Orphan drug]